PMDA approval: Evkeeza for intravenous infusion 345 mg
Applicant: Ultragenyx Japan K.K. Approval date: 2024-01-18. Type: New approval. Active ingredient(s): Evinacumab (genetical recombination).
Notes: A drug with a new active ingredient indicated for the treatment of homozygous familial hypercholesterolemia. [Orphan drug]